PMDA approval: Xyzal Tablets 5 mg
Applicant: GlaxoSmithKline K.K. Approval date: 2010-10-27. Type: New approval. Active ingredient(s): Levocetirizine hydrochloride.
Notes: A drug with a new active ingredient indicated for the treatment of allergic rhinitis, urticaria, eczema/dermatitis, pruigo and cutaneous pruritus.